PMDA approval: Talymus Ophthalmic Suspension 0.1%
Applicant: Senju Pharmaceutical Co., Ltd. Approval date: 2008-01-25. Type: New approval. Active ingredient(s): Tacrolimus hydrate.
Notes: A drug with a new route of administration indicated for the treatment of vernal conjunctivitis (in the case where anti-allergic drugs are not sufficiently effective). [Orphan drug]